PMDA approval: Scemblix Tablets 20 mg Scemblix Tablets 40 mg
Applicant: Novartis Pharma K.K. Approval date: 2022-03-28. Type: New approval. Active ingredient(s): Asciminib hydrochloride.
Notes: Drugs with a new active ingredient indicated for the treatment of chronic myelogenous leukemia with resistance or intolerance to prior drug therapies. [Orphan drug]